PMDA approval: Xarelto Tablets 2.5 mg
Applicant: Bayer Yakuhin, Ltd. Approval date: 2022-06-20. Type: New approval. Active ingredient(s): Rivaroxaban.
Notes: A drug with a new indication and a new dosage in an additional dosage form for the inhibition of thrombus/embolization formation in patients with peripheral arterial disease after lower extremity revascularization.